PMDA approval: Cylocide N Injection 400 mg Cylocide N Injection 1 g (Nippon Shinyaku Co., Ltd.) Cytarabine for I.V. Infusion 400 mg “Teva” Cytarabine for I.V. Infusion 1 g “Teva”
Applicant: Teva Takeda Pharma Ltd. Approval date: 2019-03-26. Type: Partial change. Active ingredient(s): Cytarabine.
Notes: Drugs with other characteristics indicated for the treatment prior to tumor-specific T-cell infusion therapy. [Expedited review]